PMDA approval: Irribow Tablets 2.5 μg Irribow Tablets 5 μg
Applicant: Astellas Pharma Inc. Approval date: 2008-07-16. Type: New approval. Active ingredient(s): Ramosetron hydrochloride.
Notes: Drugs with a new indication and new dosages for the treatment of diarrhea-predominant irritable bowel syndrome in men.